PMDA approval: Clomid Tablets 50 mg
Applicant: Fuji Pharma Co., Ltd. Approval date: 2022-03-11. Type: Partial change. Active ingredient(s): Clomiphene citrate.
Notes: A drug with a new indication and a new dosage for the induction of spermatogenesis in oligozoospermia. [Public knowledge-based application]